PMDA approval: Tecentriq Intravenous Infusion 1200 mg
Applicant: Chugai Pharmaceutical Co., Ltd. Approval date: 2018-01-19. Type: New approval. Active ingredient(s): Atezolizumab (genetical recombination).
Notes: A drug with a new active ingredient indicated for the treatment of unresectable advanced or recurrent non-small-cell lung cancer.